PMDA approval: Eylea Intravitreal Injection 40 mg/mL Eylea Intravitreal Injection Kit 40 mg/mL
Applicant: Bayer Yakuhin, Ltd. Approval date: 2013-11-22. Type: Partial change. Active ingredient(s): Aflibercept (genetical recombination).
Notes: Drugs with a new additional indication and a new dosage for the treatment of macular edema following central retinal vein occlusion.